PMDA approval: Savene Injectable 500 mg
Applicant: Kissei Pharmaceutical Co., Ltd. Approval date: 2014-01-17. Type: New approval. Active ingredient(s): Dexrazoxane.
Notes: A drug with a new active ingredient indicated for the treatment of anthracycline extravasation.